PMDA approval: Diagnogreen for Injection 25 mg
Applicant: Daiichi Sankyo Company, Limited. Approval date: 2023-09-25. Type: Partial change. Active ingredient(s): Indocyanine green.
Notes: A drug with a new indication and a new dosage for the visualization of extrahepatic biliary ducts. [Public knowledge-based application after preliminary assessment by the Pharmaceutical Affairs and Food Sanitation Council (PAFSC)]